PMDA approval: Rheumatrex Capsules 2 mg
Applicant: Pfizer Japan Inc. Approval date: 2019-03-26. Type: Partial change. Active ingredient(s): Methotrexate.
Notes: A drug with new additional indications and a new dosage for the treatment of plaque psoriasis in patients who have not responded sufficiently to topical therapy, or psoriatic arthritis, pustular psoriasis and erythrodermic psoriasis. [Public knowledge-based application after PAFSC's preliminary assessment]